PMDA approval: Temodal Injection 100 mg
Applicant: Schering-Plough K.K. Approval date: 2010-01-20. Type: New approval. Active ingredient(s): Temozolomide.
Notes: A drug with a new route of administration indicated for the treatment of malignant glioma.